PMDA approval: Fruzaqla Capsules 1 mg Fruzaqla Capsules 5 mg
Applicant: Takeda Pharmaceutical Company Limited. Approval date: 2024-09-24. Type: New approval. Active ingredient(s): Fruquintinib.
Notes: Drugs with a new active ingredient indicated for the treatment of unresectable advanced or recurrent colon or rectal cancer that has progressed after cancer chemotherapy.